PMDA approval: Lipiodol 480 Injection 10 mL
Applicant: Guerbet Japan K.K. Approval date: 2013-09-13. Type: Partial change. Active ingredient(s): Iodine addition products of the ethylesters of the fatty acids obtained from poppyseed oil.
Notes: A drug with a new route of administration and a new dosage for a new additional indication for the adjustment of drugs or medical devices. [Public knowledge-based application after preliminary assessment by the Pharmaceutical Affairs and Food Sanitation Council (PAFSC)]